PMDA approval: Lonsurf Combination Tablet T15 Lonsurf Combination Tablet T20
Applicant: Taiho Pharmaceutical Co., Ltd. Approval date: 2015-03-20. Type: Partial change. Active ingredient(s): (1) Trifluridine (2) Tipiracil hydrochloride.
Notes: Drugs with a revised indication for the treatment of unresectable advanced or recurrent colorectal cancer. [Expedited review]